PMDA approval: Isturisa Tablets 1 mg Isturisa Tablets 5 mg
Applicant: Recordati Rare Diseases Japan K.K. Approval date: 2021-03-23. Type: New approval. Active ingredient(s): Osilodrostat phosphate.
Notes: Drugs with a new active ingredient indicated for the treatment of Cushing's syndrome (when surgical therapies are not sufficiently effective or are difficult to be performed).